PMDA approval: Infliximab BS for I.V. Infusion 100 mg “Nichiiko” (Nichi-Iko Pharmaceutical Co., Ltd.) Infliximab BS for I.V. Infusion 100 mg “AYUMI”
Applicant: Yakuhan Pharmaceutical Co., Ltd. Approval date: 2017-09-27. Type: New approval. Active ingredient(s): Infliximab (genetical recombination) [Infliximab biosimilar 2].
Notes: Follow-on biologics indicated for the treatment of rheumatoid arthritis, psoriasis, Crohn’s disease, and ulcerative colitis.